PMDA approval: Mektovi Tablets 15 mg
Applicant: Ono Pharmaceutical Co., Ltd. Approval date: 2019-01-08. Type: New approval. Active ingredient(s): Binimetinib.
Notes: A drug with a new active ingredient indicated for the treatment of unresectable melanoma with BRAF gene mutation. [Orphan drug]